PMDA approval: Kesimpta for s.c.injection 20 mg Pen
Applicant: Novartis Pharma K.K. Approval date: 2021-03-23. Type: New approval. Active ingredient(s): Ofatumumab (genetical recombination).
Notes: A drug with a new route of administration and new indications for the prevention of relapse and for delaying the accumulation of physical disability in relapsing-remitting multiple sclerosis (RRMS) and active secondary progressive multiple sclerosis (active SPMS). [Orphan drug]